PMDA approval: Repatha SC Injection 140 mg Syringe Repatha SC Injection 140 mg Pen Repatha SC Injection 420 mg Auto Mini-doser
Applicant: Amgen Astellas BioPharma K.K. Approval date: 2019-06-18. Type: Partial change. Active ingredient(s): Evolocumab (genetical recombination).
Notes: Drugs with a new additional indication for the treatment of familial hypercholesterolemia and hypercholesterolemia (for use only in patients who are at higher risk of developing cardiovascular events, and in whom treatment with HMG-CoA reductase inhibitors is not suitable).